PMDA approval: Arexvy Intramuscular Injection
Applicant: GlaxoSmithKline K.K. Approval date: 2024-11-22. Type: Partial change. Active ingredient(s): Recombinant respiratory syncytial virus PreF3 antigen.
Notes: A drug with a new dosage indicated for the prevention of disease caused by RS virus infection in individuals aged 50 years or older who are considered to be at a high risk of developing severe RS virus infection.